PMDA approval: Tracleer Tablets 62.5 mg
Applicant: Actelion Pharmaceuticals Japan Ltd. Approval date: 2012-11-21. Type: Partial change. Active ingredient(s): Bosentan hydrate.
Notes: A drug with a new additional indication for the treatment of pulmonary arterial hypertension (WHO functional class II). [Orphan drug]